PMDA approval: AtvagoReverse Intravenous Injection Syringe 3 mL AtvagoReverse Intravenous Injection Syringe 6 mL
Applicant: Terumo Corporation. Approval date: 2008-07-16. Type: New approval. Active ingredient(s): Neostigmine methylsulfate/ atropine sulfate hydrate.
Notes: New combination drugs indicated for reversal of nondepolarizing muscle relaxants.